PMDA approval: Taxol Injection 30 mg Taxol Injection 100 mg
Applicant: Bristol-Myers K.K. Approval date: 2007-12-12. Type: Partial change. Active ingredient(s): Paclitaxel.
Notes: Drugs with a new dosage indicated for the treatment of breast cancer, with once-weekly dosing continuously for 6 weeks followed by a 2-week withdrawal period.